PMDA approval: Romiplate for S.C. Injection 250 µg
Applicant: Kyowa Hakko Kirin Co., Ltd. Approval date: 2019-06-18. Type: Partial change. Active ingredient(s): Romiplostim (genetical recombination).
Notes: A drug with a new indication and a new dosage for the treatment of aplastic anemia in patients who have not responded sufficiently to conventional treatments.